PMDA approval: Erwinase for Intramuscular Injection 10000
Applicant: Ohara Pharmaceutical Co., Ltd. Approval date: 2016-12-19. Type: New approval. Active ingredient(s): Crisantaspase.
Notes: A drug with a new active ingredient indicated for the treatment of acute leukemia (including blast crisis of chronic leukemia) and malignant lymphoma (only for patients who experienced hypersensitivity to L-asparaginase preparations).